PMDA approval: Ditripentat-Cal Injection 1000 mg Zinc-Tripentat Injection 1055 mg
Applicant: Nihon Medi-Physics Co., Ltd. Approval date: 2011-07-01. Type: New approval. Active ingredient(s): Pentetate calcium trisodium Pentetate zinc trisodium.
Notes: Drugs with a new active ingredient indicated for the reduction of internal contamination with transuranium elements (plutonium, americium, and curium). [Expedited review]